PMDA approval: Betanis Tablet 25 mg Betanis Tablet 50 mg
Applicant: Astellas Pharma Inc. Approval date: 2011-07-01. Type: New approval. Active ingredient(s): Mirabegron.
Notes: Drugs with a new active ingredient indicated for the treatment of urinary urgency, urinary frequency, and urge urinary incontinence associated with overactive bladder.